PMDA approval: Cylocide Injection 20 mg Cylocide Injection 40 mg Cylocide Injection 60 mg Cylocide Injection 100 mg Cylocide Injection 200 mg
Applicant: Nippon Shinyaku Co., Ltd. Approval date: 2020-08-21. Type: Partial change. Active ingredient(s): Cytarabine.
Notes: Drugs with a new dosage indicated for the treatment of acute leukemia (including erythroleukemia and blast crisis of chronic myeloid leukemia). [Public knowledge-based application]